PMDA approval: Ronapreve for Intravenous Infusion Set 300 Ronapreve for Intravenous Infusion Set 1332
Applicant: Chugai Pharmaceutical Co., Ltd. Approval date: 2021-07-19. Type: New approval. Active ingredient(s): Casirivimab (genetical recombination), Imdevimab (genetical recombination).
Notes: Drugs with new active ingredients indicated for the treatment of disease caused by SARS-CoV-2 infection (COVID-19). [Drug pertaining to Special Approval for Emergency]